PMDA approval: Paclitaxel Inj. 30 mg/5 mL "NK" Paclitaxel Inj. 100 mg/16.7 mL "NK"
Applicant: Nippon Kayaku Co., Ltd. Approval date: 2022-09-26. Type: Partial change. Active ingredient(s): Paclitaxel.
Notes: Drugs with a new dosage indicated for thetreatment of advanced or recurrent cervical cancer. [Public knowledge-based application]